PMDA approval: Yuvanci Combination Tablets
Applicant: Janssen Pharmaceutical K.K. Approval date: 2024-09-24. Type: New approval. Active ingredient(s): Macitentan/Tadalafil.
Notes: A new combination drug indicated for the treatment of pulmonary arterial hypertension.